PMDA approval: Cellcept Capsules 250 Cellcept Powder for Oral Suspension 31.8%
Applicant: Chugai Pharmaceutical Co., Ltd. Approval date: 2024-06-24. Type: Partial change. Active ingredient(s): Mycophenolate mofetil.
Notes: Drugs with a new indication and a new dosage for the treatment of systemic sclerosis-associated interstitial lung disease. [Public knowledge-based application after PAFSC's preliminary assessment]